PMDA approval: Laserphyrin 100 mg for Injection
Applicant: Meiji Seika Pharma Co., Ltd. Approval date: 2013-09-20. Type: Partial change. Active ingredient(s): Talaporfin sodium.
Notes: A drug with a new additional indication and a new dosage for the treatment of primary malignant brain tumor (only for the case where surgical excision of tumor is performed). [Orphan drug]